PMDA approval: Jivi for iv injection 250 Jivi for iv injection 500 Jivi for iv injection 1000 Jivi for iv injection 2000 Jivi for iv injection 3000
Applicant: Bayer Yakuhin, Ltd. Approval date: 2018-09-21. Type: New approval. Active ingredient(s): Damoctocog alfa pegol (genetical recombination).
Notes: Drugs with a new active ingredient indicated for the control of bleeding tendency in patients with blood coagulation factor VIII deficiency.